PMDA approval: Allermist 27.5 μg 56 metered Nasal Spray
Applicant: GlaxoSmithKline K.K. Approval date: 2009-04-22. Type: New approval. Active ingredient(s): Fluticasone furoate.
Notes: A drug with a new active ingredient indicated for the treatment of allergic rhinitis.